PMDA approval: Keytruda Injection 20 mg Keytruda Injection 100 mg
Applicant: MSD K.K. Approval date: 2017-12-25. Type: Partial change. Active ingredient(s): Pembrolizumab (genetical recombination).
Notes: Drugs with a new additional indication for the treatment of unresectable urothelial cancer which progressed after cancer chemotherapy. [Priority review]